PMDA approval: Nailin Capsules 100 mg
Applicant: Sato Pharmaceutical Co., Ltd. Approval date: 2018-01-19. Type: New approval. Active ingredient(s): Fosravuconazole L-lysine ethanolate.
Notes: A drug with a new active ingredient indicated for the treatment of onychomycosis due to Trichophyton species.